PMDA approval: Penicillin G Potassium 200000 Units for Injection Penicillin G Potassium 1000000 Units for Injection
Applicant: Meiji Seika Pharma Co., Ltd. Approval date: 2012-02-22. Type: Partial change. Active ingredient(s): Benzylpenicillin potassium.
Notes: Drugs with a new route of administration and a new dosage for a new additional indication for the treatment of syphilis. [Public knowledge-based application after PAFSC's preliminary assessment]